PMDA approval: Regpara Tablets 25 mg Regpara Tablets 75 mg
Applicant: Kirin Pharma Co., Ltd. Approval date: 2007-10-19. Type: New approval. Active ingredient(s): Cinacalcet hydrochloride.
Notes: A drug containing a new active ingredient indicated for the treatment of secondary hyperparathyroidism in patients on maintenance dialysis.